PMDA approval: Cuvitru 20% S.C. Injection 1 g/5 mL Cuvitru 20% S.C. Injection 2 g/10 mL Cuvitru 20% S.C. Injection 4 g/20 mL Cuvitru 20% S.C. Injection 8 g/40 mL Cuvitru 20% S.C. Injection 10 g/50 mL
Applicant: Takeda Pharmaceutical Company, Limited. Approval date: 2023-09-25. Type: New approval. Active ingredient(s): Human normal immunoglobulin G (pH4-treated acidic normal human immunoglobulin [subcutaneous injection]).
Notes: Drugs with a new active ingredient indicated for the treatment of agammaglobulinemia or hypogammaglobulinemia.